PMDA approval: Orkedia Tablets 1 mg Orkedia Tablets 2 mg
Applicant: Kyowa Kirin Co., Ltd. Approval date: 2019-12-20. Type: Partial change. Active ingredient(s): Evocalcet.
Notes: Drugs with new additional indications and a new dosage for the treatment of hypercalcemia in patients with parathyroid carcinoma or patients with primary hyperparathyroidism who are unable to undergo parathyroidectomy or relapse after parathyroidectomy. [Orphan drug]